PMDA approval: Tribik
Applicant: The Research Foundation for Microbial Diseases of Osaka University. Approval date: 2016-02-29. Type: Partial change. Active ingredient(s): Adsorbed diphtheria-purified pertussis-tetanus combined vaccine.
Notes: A drug with a new dosage indicated for the prevention of pertussis, diphtheria, and tetanus.